PMDA approval: Deberza Tablets 20 mg (Kowa Company, Ltd.) Apleway 20 mg Tablets
Applicant: Sanofi K.K. Approval date: 2014-03-24. Type: New approval. Active ingredient(s): Tofogliflozin hydrate.
Notes: Drugs with a new active ingredient indicated for the treatment of type 2 diabetes mellitus.